PMDA approval: Efluelda intramuscular injection
Applicant: Sanofi K.K. Approval date: 2024-12-27. Type: New approval. Active ingredient(s): Influenza virus (type A and type B) HA fraction.
Notes: A drug with a new active ingredient indicated for the prevention of influenza.